PMDA approval: Inrebic Capsules 100 mg
Applicant: Bristol-Myers Squibb K.K. Approval date: 2025-06-24. Type: New approval. Active ingredient(s): Fedratinib hydrochloride hydrate.
Notes: A drug with a new active ingredient indicated for the treatment of myelofibrosis. [Orphan drug]